PMDA approval: Rozlytrek Capsules 100 mg Rozlytrek Capsules 200 mg
Applicant: Chugai Pharmaceutical Co., Ltd. Approval date: 2020-02-21. Type: Partial change. Active ingredient(s): Entrectinib.
Notes: Drugs with a new indication and a new dosage for the treatment of ROS1 fusion gene-positive unresectable advanced or recurrent non-small-cell lung cancer.